PMDA approval: Cymbalta Capsules 20 mg Cymbalta Capsules 30 mg
Applicant: Shionogi & Co., Ltd. Approval date: 2016-12-19. Type: Partial change. Active ingredient(s): Duloxetine hydrochloride.
Notes: Drugs with a new additional indication for the treatment of pain associated with osteoarthritis.